PMDA approval: Norditropin S Injection 5 mg Norditropin S Injection 10 mg Norditropin NordiFlex Injection 5 mg Norditropin NordiFlex Injection 10 mg Norditropin NordiFlex Injection 15 mg
Applicant: Novo Nordisk Pharma Ltd. Approval date: 2009-04-22. Type: Partial change. Active ingredient(s): Somatropin (genetical recombination).
Notes: Drugs with a new additional indication and a new dosage for the treatment of adult growth hormone deficiency (for use only in severe cases).